PMDA approval: Intuniv Tablets 1 mg Intuniv Tablets 3 mg
Applicant: Shionogi & Co., Ltd. Approval date: 2017-03-30. Type: New approval. Active ingredient(s): Guanfacine hydrochloride.
Notes: Drugs with a new active ingredient indicated for the treatment of pediatric attention deficit/hyperactivity disorder (AD/HD).